PMDA approval: (1) Pegfilgrastim BS Subcutaneous Injection 3.6 mg "Mochida" (2) Pegfilgrastim BS Subcutaneous Injection 3.6 mg "Nipro"
Applicant: [1] Mochida Pharmaceutical Co., Ltd. [2] Mochida Pharmaceutical Sales Co., Ltd. Approval date: 2023-09-25. Type: New approval. Active ingredient(s): Pegfilgrastim (genetical recombination) [pegfilgrastim biosimilar 1].
Notes: Biosimilars indicated for the prevention of febrile neutropenia in patients receiving cancer chemotherapy.